PMDA approval: Unitalc Intrapleural 4 g
Applicant: Nobelpharma Co., Ltd. Approval date: 2013-09-20. Type: New approval. Active ingredient(s): Sterile talc.
Notes: A drug with a new active ingredient indicated for the suppression of recurrence of malignant pleural effusions.